PMDA approval: Rasilez Tablets 150 mg
Applicant: Novartis Pharma K.K. Approval date: 2009-07-07. Type: New approval. Active ingredient(s): Aliskiren fumarate.
Notes: A drug with a new active ingredient indicated for the treatment of hypertension.